PMDA approval: Gardasil Aqueous Suspension for Intramuscular Injection Gardasil Aqueous Suspension for Intramuscular Injection Syringe
Applicant: MSD K.K. Approval date: 2011-07-01. Type: New approval. Active ingredient(s): Recombinant adsorbed quadrivalent human papillomavirus virus-like particle vaccine (yeast origin).
Notes: Drugs with a new active ingredient indicated for the prevention of cervical cancer and its precursor lesions, vulvar intraepithelial neoplasia, vaginal intraepithelial neoplasia, and condyloma acuminatum caused by infection with human papillomavirus types 6, 11, 16 and 18.